PMDA approval: Xolair for S.C. Injection 75 mg Xolair for S.C. Injection 150 mg Xolair for S.C. Injection 75 mg Syringe Xolair for S.C. Injection 150 mg Syringe
Applicant: Novartis Pharma K.K. Approval date: 2019-12-11. Type: Partial change. Active ingredient(s): Omalizumab (genetical recombination).
Notes: Drugs with a new additional indication and a new dosage for the treatment of seasonal allergic rhinitis (for use only in severe or the most severe patients who have not responded sufficiently to conventional therapies).